PMDA approval: Orencia for I.V. Infusion 250 mg
Applicant: Bristol-Myers Squibb K.K. Approval date: 2018-02-23. Type: Partial change. Active ingredient(s): Abatacept (genetical recombination).
Notes: A drug with a new additional indication and a new dosage indicated for the treatment of polyarticular-course juvenile idiopathic arthritis in patients who have not responded sufficiently to conventional therapies.